PMDA approval: Zerbaxa Combination for Intravenous Drip Infusion
Applicant: MSD K.K. Approval date: 2019-12-20. Type: Partial change. Active ingredient(s): Ceftolozane sulfate/Tazobactam sodium.
Notes: A drug with new additional indications and a new dosage for the treatment of sepsis and pneumonia caused by Serratia and Haemophilus influenzae.